PMDA approval: Aricept Tablets 3 mg Aricept Tablets 5 mg Aricept Tablets 10 mg Aricept D Tablets 3 mg Aricept D Tablets 5 mg Aricept D Tablets 10 mg Aricept Fine Granules 0.5%
Applicant: Eisai Co., Ltd. Approval date: 2007-08-23. Type: Partial change. Active ingredient(s): Donepezil hydrochloride.
Notes: Drugs with a new indication and dosage for patients with severe dementia of the Alzheimer’ s type.